PMDA approval: Naruvein Injection 2 mg Naruvein Injection 20 mg
Applicant: Daiichi Sankyo Propharma Co., Ltd. Approval date: 2018-01-19. Type: New approval. Active ingredient(s): Hydromorphone hydrochloride.
Notes: Drugs with a new route of administration indicated for management of moderate to severe pain in various types of cancer.